PMDA approval: Ozex Fine Granules 15% for Pediatric
Applicant: Toyama Chemical Co., Ltd. Approval date: 2017-03-02. Type: Partial change. Active ingredient(s): Tosufloxacin tosilate hydrate.
Notes: A drug with a new additional indication for the treatment of mycoplasma pneumonia caused by Mycoplasma pneumoniae.